PMDA approval: MaQaid Intravitreal Injection 40 mg
Applicant: Wakamoto Co., Ltd. Approval date: 2010-10-27. Type: New approval. Active ingredient(s): Triamcinolone acetonide.
Notes: A drug with a new route of administration indicated for the visualization of the vitreous body during vitreous surgery.